PMDA approval: Sarclisa 100 mg I.V. Infusion Sarclisa 500 mg I.V. Infusion
Applicant: Sanofi K.K. Approval date: 2021-11-25. Type: Partial change. Active ingredient(s): Isatuximab (genetical recombination).
Notes: Drugs with a new dosage indicated for the treatment of relapsed or refractory multiple myeloma.